PMDA approval: Joyclu 30 mg Intra-articular Injection
Applicant: Seikagaku Corporation. Approval date: 2021-03-23. Type: New approval. Active ingredient(s): Diclofenac etalhyaluronate sodium.
Notes: A drug with a new active ingredient indicated for the treatment of knee and hip osteoarthritis.